PMDA approval: Bydureon for Subcutaneous Injection 2 mg
Applicant: Eli Lilly Japan K.K. Approval date: 2012-03-30. Type: New approval. Active ingredient(s): Exenatide.
Notes: A drug with a new additional indication and a new dosage in a new dosage form for the treatment of type 2 diabetes mellitus.